PMDA approval: Lagnos NF Jelly for Oral Administration Divided Pack 12 g
Applicant: Sanwa Kagaku Kenkyusho Co., Ltd. Approval date: 2018-09-21. Type: New approval. Active ingredient(s): Lactulose crystal.
Notes: A drug with a new additional indication and a new dosage in an additional dosage form indicated for the treatment of chronic constipation (excluding constipation due to organic diseases).